PMDA approval: Comirnaty Intramuscular Injection for 6 months to 4 years old
Applicant: Pfizer Japan Inc. Approval date: 2022-10-05. Type: New approval. Active ingredient(s): Tozinameran.
Notes: A drug with a new additional pediatric dosage in an additional dosage form indicated for the prevention of disease caused by SARS-CoV-2 infection (COVID-19). [Drug pertaining to Special Approval for Emergency]